PMDA approval: Spinraza Intrathecal injection 12 mg
Applicant: Biogen Japan Ltd. Approval date: 2017-09-22. Type: Partial change. Active ingredient(s): Nusinersen sodium.
Notes: A drug with a revised indication and a new dosage for the treatment of spinal muscular atrophy. [Orphan drug]